PMDA approval: Opdivo Intravenous Infusion 20 mg Opdivo Intravenous Infusion 100 mg Opdivo Intravenous Infusion 120 mg Opdivo Intravenous Infusion 240 mg
Applicant: Ono Pharmaceutical Co., Ltd. Approval date: 2021-11-25. Type: Partial change. Active ingredient(s): Nivolumab (genetical recombination).
Notes: 1) Drugs with a new indication and a new dosage for the treatment of unresectable advanced or recurrent gastric cancer. 2) Drugs with a new indication and a new dosage for postoperative adjuvant therapy for esophageal cancer.